PMDA approval: Malarone Combination Tablets Malarone Pediatric Combination Tablets
Applicant: GlaxoSmithKline K.K. Approval date: 2016-03-28. Type: Partial change. Active ingredient(s): Atovaquone / Proguanil hydrochloride.
Notes: A drug with a revised pediatric dosage and an additional dosage form indicated for the treatment and prevention of malaria.